PMDA approval: Endoxan Tablets 50 mg
Applicant: Shionogi & Co., Ltd. Approval date: 2011-09-16. Type: Partial change. Active ingredient(s): Cyclophosphamide hydrate.
Notes: A drug with a new additional indication and a new dosage for the treatment of nephrotic syndrome (for use only in patients who have not sufficiently responded to adequate treatment with corticosteroids). [Public knowledge-based application after PAFSC's preliminary assessment]